PMDA approval: Propess Vaginal Inserts 10 mg
Applicant: Ferring Pharmaceuticals Co., Ltd. Approval date: 2020-01-23. Type: New approval. Active ingredient(s): Dinoprostone.
Notes: A drug with a new route of administration indicated for the initiation of cervical ripening in patients with unfavourable cervix from 37 completed weeks of gestation.